PMDA approval: Carbostar 2 · L for Dialysis Carbostar 2 · P for Dialysis
Applicant: Yoshindo Inc. Approval date: 2025-03-27. Type: New approval. Active ingredient(s): N/A for this combination drug.
Notes: Combination prescription drugs with similar formulations to be used as perfusion fluids for hemodialysis in patients with chronic renal failure (use in any of the following cases: • When the patient’s blood sugar level is not controlled well with sugarless dialysate. • When dialysate with a high calcium concentration may cause hypercalcemia).